PMDA approval: Keytruda Injection 100 mg
Applicant: MSD K.K. Approval date: 2024-09-24. Type: Partial change. Active ingredient(s): Pembrolizumab (genetical recombination).
Notes: A drug with a new indication for the treatment of unresectable urothelial carcinoma. [Priority review]